PMDA approval: Actemra 80 mg for Intravenous Infusion Actemra 200 mg for Intravenous Infusion Actemra 400 mg for Intravenous Infusion
Applicant: Chugai Pharmaceutical Co., Ltd. Approval date: 2022-01-21. Type: Partial change. Active ingredient(s): Tocilizumab (genetical recombination).
Notes: Drugs with a new indication and a new dosage for the treatment of pneumonia caused by SARS-CoV-2 (only for the patients requiring supplemental oxygen). [Priority review]